PMDA approval: Takepron Capsules 15 Takepron OD Tablets 15
Applicant: Takeda Pharmaceutical Company Limited. Approval date: 2010-07-23. Type: Partial change. Active ingredient(s): Lansoprazole.
Notes: Drugs with a new additional indication and a new dosage for prevention of recurrence of gastric ulcer or duodenal ulcer in patients treated with low-dose aspirin.